PMDA approval: Keytruda Injection 100 mg
Applicant: MSD K.K. Approval date: 2024-08-28. Type: Partial change. Active ingredient(s): Pembrolizumab (genetical recombination).
Notes: A drug with a new indication and a new dosage for the preoperative and postoperative adjuvant treatment for non-small cell lung cancer.